PMDA approval: Azilect Tablets 0.5 mg Azilect Tablets 1 mg
Applicant: Takeda Pharmaceutical Company Limited. Approval date: 2018-03-23. Type: New approval. Active ingredient(s): Rasagiline mesilate.
Notes: Drugs with a new active ingredient indicated for the treatment of Parkinson’s disease.